PMDA approval: Rabipur Intramuscular Injection
Applicant: GlaxoSmithKline K.K. Approval date: 2019-03-26. Type: New approval. Active ingredient(s): Freeze-dried inactivated tissue culture rabies vaccine.
Notes: A drug with a new active ingredient indicated for the pre-exposure and post-exposure prophylaxis against rabies.